PMDA approval: Jemina Tablets
Applicant: Nobelpharma Co., Ltd. Approval date: 2018-07-02. Type: New approval. Active ingredient(s): Levonorgestrel/Ethinylestradiol.
Notes: A new combination drug indicated for the treatment of dysmenorrhea.